PMDA approval: Taltz 80 mg Syringe for SC Injection Taltz 80 mg Auto-Injector for SC Injection
Applicant: Eli Lilly Japan K.K. Approval date: 2018-08-21. Type: Partial change. Active ingredient(s): Ixekizumab (genetical recombination).
Notes: Drugs with a new dosage indicated for the treatment of plaque psoriasis, psoriatic arthritis, pustular psoriasis, and erythrodermic psoriasis in patients who have not responded sufficiently to conventional therapies.